PMDA approval: AmBisome 50 mg for Intravenous Drip Infusion
Applicant: Dainippon Sumitomo Pharma Co., Ltd. Approval date: 2009-06-17. Type: Partial change. Active ingredient(s): Amphotericin B.
Notes: A drug with new additional indications and a new dosage for the treatment of fungal infections caused by Mucor species, Absidia species, Rhizopus species, Rhizomucor species, Cladosporium species, Cladophialophora species, Fonsecaea species, Phialophora species, Exophiala species, Coccidioides species, Histoplasma species, and Blastomyces species and visceral leishmaniasis.